PMDA approval: Carboplatin Injection 50 mg "NK" Carboplatin Injection 150 mg "NK" Carboplatin Injection 450 mg "NK"
Applicant: Mylan Seiyaku Ltd. Approval date: 2021-08-25. Type: Partial change. Active ingredient(s): Carboplatin.
Notes: Drugs with a new dosage indicated for the treatment of breast cancer.